PMDA approval: Bup-4 Tablet 10 Bup-4 Tablet 20 Bup-4 Fine Granule 2%
Applicant: Taiho Pharmaceutical Co., Ltd. Approval date: 2009-12-18. Type: Partial change. Active ingredient(s): Propiverine hydrochloride.
Notes: Drugs with a new additional indication for the treatment of urgency of urination, pollakiuria, and urge urinary incontinence associated with overactive bladder.